PMDA approval: ABILIFY Prolonged Release Aqueous Suspension for IM Injection 300 mg ABILIFY Prolonged Release Aqueous Suspension for IM Injection 400 mg ABILIFY Prolonged Release Aqueous Suspension for IM Injection 300 mg Syringe ABILIFY Prolonged Release Aqueous Suspension for IM Injection 400 mg Syringe
Applicant: Otsuka Pharmaceutical Co., Ltd. Approval date: 2015-03-26. Type: New approval. Active ingredient(s): Aripiprazole hydrate.
Notes: Drugs with a new route of administration indicated for the treatment of schizophrenia.